PMDA approval: Treakisym Injection 100 mg
Applicant: SymBio Pharmaceuticals Limited. Approval date: 2016-08-26. Type: Partial change. Active ingredient(s): Bendamustine hydrochloride.
Notes: A drug with a new additional indication and a new dosage for the treatment of chronic lymphocytic leukemia. [Orphan drug]